PMDA approval: Humira 40 mg for S.C. Injection Syringe 0.4 mL Humira 80 mg for S.C. Injection Syringe 0.8 mL Humira 40 mg for S.C. Injection in pre-filled pen 0.4 mL Humira 80 mg for S.C. Injection in pre-filled pen 0.8 mL
Applicant: AbbVie GK. Approval date: 2020-05-29. Type: Partial change. Active ingredient(s): Adalimumab (genetical recombination).
Notes: Drugs with a new dosage indicated for the treatment of hidradenitis suppurativa. [Orphan drug]